PMDA approval: Novastan HI Inj. 10 mg/2mL (Mitsubishi Tanabe Pharma Corporation) Slonnon HI Injection 10 mg/2mL
Applicant: Daiichi Sankyo Co., Ltd. Approval date: 2008-07-16. Type: Partial change. Active ingredient(s): Argatroban hydrate.
Notes: Drugs with a new indication and a new dosage for prophylaxis of thrombosis in patients with heparin-induced thrombocytopenia (HIT) type II. [Orphan drug]